PMDA approval: Ebglyss Subcutaneous Injection 250 mg Syringes Ebglyss Subcutaneous Injection 250 mg Autoinjectors
Applicant: Eli Lilly Japan K.K. Approval date: 2024-01-18. Type: New approval. Active ingredient(s): Lebrikizumab (genetical recombination).
Notes: Drugs with a new active ingredient indicated for the treatment of atopic dermatitis in patients who have not responded sufficiently to conventional treatments.